PMDA approval: Yorvipath Subcutaneous Injection 168 µg Pen Yorvipath Subcutaneous Injection 294 µg Pen Yorvipath Subcutaneous Injection 420 µg Pen
Applicant: Teijin Pharma Limited. Approval date: 2025-08-25. Type: New approval. Active ingredient(s): Palopegteriparatide.
Notes: Drugs with a new active ingredient indicated for the treatment of hypoparathyroidism. [Orphan drug]